PMDA approval: Immucyst Intravesical 81 mg
Applicant: Nippon Kayaku Co., Ltd. Approval date: 2010-08-20. Type: Partial change. Active ingredient(s): Freeze-dried bacillus of Calmette and Guerin for intravesical use (Connaught strain).
Notes: A drug with a new additional dosage indicated for the treatment of superficial bladder cancer and carcinoma in situ of bladder.